PMDA approval: Rezaltas Combination Tablets LD Rezaltas Combination Tablets HD
Applicant: Daiichi Sankyo Co., Ltd. Approval date: 2010-01-20. Type: New approval. Active ingredient(s): Olmesartan medoxomil/ azelnidipine.
Notes: New combination drugs indicated for the treatment of hypertension.